PMDA approval: Cimzia 200 mg Syringe for S.C. Injection
Applicant: UCB Japan Co. Ltd. Approval date: 2015-05-26. Type: Partial change. Active ingredient(s): Certolizumab pegol (genetical recombination).
Notes: A drug with a revised indication for the treatment of rheumatoid arthritis (including prevention of structural joint damage).